PMDA approval: Luconac Solution 5 ％
Applicant: Sato Pharmaceutical Co., Ltd. Approval date: 2016-01-22. Type: New approval. Active ingredient(s): Luliconazole.
Notes: A drug with a new indication in a new dosage form indicated for the treatment of tinea unguium.